PMDA approval: Stribild Combination Tab.
Applicant: Japan Tobacco Inc. Approval date: 2013-03-25. Type: New approval. Active ingredient(s): Elvitegravir/cobicistat/ emtricitabine/tenofovir disoproxil fumarate.
Notes: A new combination drug with a new active ingredient indicated for the treatment of HIV-1 infection. [Orphan drug]